PMDA approval: Humira 40mg for S.C. Injection
Applicant: Abbott Japan Co., Ltd. Approval date: 2008-04-16. Type: New approval. Active ingredient(s): Adalimumab (genetical recombination).
Notes: A drug containing a new active ingredient indicated for the treatment of rheumatoid arthritis (for use only when conventional therapies are not sufficiently effective).